PMDA approval: (1) Brilinta Tablets 60 mg (2) Brilinta Tablets 90 mg
Applicant: AstraZeneca K.K. Approval date: 2016-09-28. Type: New approval. Active ingredient(s): Ticagrelor.
Notes: Drugs with a new active ingredient indicated for the treatment of: (1) Old myocardial infarction at especially high risk of developing atherothrombosis with at least one of the following risk factors: age of 65 years or older, with diabetes mellitus requiring drug therapy, history of two or more episodes of myocardial infarction, angiography-confirmed multivessel coronary artery disease, or non-end-stage chronic renal dysfunction. (2) Acute coronary syndrome (unstable angina, non-ST-segment elevation myocardial infarction, and ST-segment elevation myocardial infarction) for which percutaneous coronary intervention (PCI) is indicated. (provided that dual antiplatelet therapy including aspirin is appropriate but the administration of other antiplatelet drugs in combination with aspirin is not suitable for the patient.)